PMDA approval: Kyprolis for Intravenous Injection 10 mg Kyprolis for Intravenous Injection 40 mg
Applicant: Ono Pharmaceutical Co., Ltd. Approval date: 2019-11-22. Type: Partial change. Active ingredient(s): Carfilzomib.
Notes: Drugs with a new dosage indicated for the treatment of relapsed or refractory multiple myeloma. [Orphan drug]